PMDA approval: Imdelltra for I.V. Infusion 1 mg Imdelltra for I.V. Infusion 10 mg
Applicant: Amgen K.K. Approval date: 2024-12-27. Type: New approval. Active ingredient(s): Tarlatamab (genetical recombination).
Notes: Drugs with a new active ingredient indicated for the treatment of small cell lung cancer that has progressed after cancer chemotherapy. [Orphan drug]